PMDA approval: Etanercept BS 10 mg for S.C. Inj. "MA" Etanercept BS 25 mg for S.C. Inj. "MA" Etanercept BS 25 mg Syringe 0.5 mL for S.C. Inj. "MA" Etanercept BS 50 mg Syringe 1.0 mL for S.C. Inj. "MA" Etanercept BS 50 mg PEN 1.0 mL for S.C. Inj. "MA"
Applicant: Mochida Pharmaceutical Co., Ltd. Approval date: 2018-01-19. Type: New approval. Active ingredient(s): Etanercept (genetical recombination) [etanercept biosimilar 1].
Notes: Follow-on biologics indicated for the treatment of rheumatoid arthritis (including the prevention of structural joint damage) and polyarticular-course juvenile idiopathic arthritis in patients who have not responded sufficiently to conventional therapies.